PMDA approval: Rasuritek 1.5 mg for I.V. Infusion Rasuritek 7.5 mg for I.V. Infusion
Applicant: Sanofi-Aventis K.K. Approval date: 2009-10-16. Type: New approval. Active ingredient(s): Rasburicase (genetical recombination).
Notes: Drugs with a new active ingredient indicated for the treatment of hyperuricemia in patients receiving cancer chemotherapy.